PMDA approval: Epoprostenol ACT 0.5 mg Epoprostenol ACT 1.5 mg
Applicant: Actelion Pharmaceuticals Japan Ltd. Approval date: 2017-03-02. Type: Partial change. Active ingredient(s): Epoprostenol sodium.
Notes: Drugs with a new additional pediatric dosage indicated for the treatment of pulmonary arterial hypertension.